PMDA approval: Synagis 50 mg solution for Intramuscular Administration Synagis 100 mg solution for Intramuscular Administration
Applicant: AstraZeneca K.K. Approval date: 2024-03-26. Type: Partial change. Active ingredient(s): Palivizumab (genetical recombination).
Notes: Drugs with a new indication for the prevention of serious lower respiratory tract disease caused by respiratory syncytial (RS) virus infection in neonates, infants, and children aged 24 months and younger with pulmonary hypoplasia, airway stenosis, congenital esophageal atresia, congenital metabolic disorder, or neuromuscular disease at the early stage of the RS virus season. [Priority review]